PMDA approval: Equfina Tablets 50 mg
Applicant: Meiji Seika Pharma Co., Ltd. Approval date: 2019-09-20. Type: New approval. Active ingredient(s): Safinamide mesilate.
Notes: A drug with a new active ingredient indicated for the improvement of wearing-off phenomenon in patients with Parkinson’s disease under the treatment with levodopa-containing preparations.